PMDA approval: Campto 40 mg for I.V. Infusion Campto 100 mg for I.V. Infusion
Applicant: Yakult Honsha Co., Ltd. Approval date: 2013-12-20. Type: Partial change. Active ingredient(s): Irinotecan hydrochloride hydrate.
Notes: Drugs with a new additional indication and a new dosage for the treatment of unresectable pancreatic cancer. [Priority review], [Expedited review]